PMDA approval: Lamictal Tablets 2 mg for Children Lamictal Tablets 5 mg for Children Lamictal Tablets 25 mg Lamictal Tablets 100 mg
Applicant: GlaxoSmithKline K.K. Approval date: 2015-09-24. Type: Partial change. Active ingredient(s): Lamotrigine.
Notes: Drugs with a new additional indication and a new dosage for use in the monotherapy for the treatment of typical absence seizures in patients with epilepsy.